PMDA approval: Glufast Tablets 5 mg Glufast Tablets 10 mg
Applicant: Kissei Pharmaceutical Co., Ltd. Approval date: 2007-05-24. Type: Partial change. Active ingredient(s): Mitiglinide calcium hydrate.
Notes: Drugs with a new indication for use in combination with α-glucosidase inhibitors to reduce glycemic excursions in patients with type 2 diabetes mellitus in whom diet and exercise therapy is not sufficiently effective.